PMDA approval: Revolade Tablets 12.5 mg Revolade Tablets 25 mg
Applicant: GlaxoSmithKline K.K. Approval date: 2010-10-27. Type: New approval. Active ingredient(s): Eltrombopag olamine.
Notes: Drugs with a new active ingredient indicated for the treatment of chronic idiopathic thrombocytopenic purpura. [Orphan drug]